PMDA approval: J Zoloft Tablets 25 mg J Zoloft Tablets 50 mg J Zoloft Tablets 100 mg J Zoloft OD Tablets 25 mg J Zoloft OD Tablets 50 mg J Zoloft OD Tablets 100 mg
Applicant: Pfizer Japan Inc. Approval date: 2015-03-20. Type: Partial change. Active ingredient(s): Sertraline hydrochloride.
Notes: Drugs with a new additional indication for the treatment of posttraumatic stress disorder.